PMDA approval: Livazebe Combination Tablets LD Livazebe Combination Tablets HD
Applicant: Kowa Company, Ltd. Approval date: 2022-09-26. Type: New approval. Active ingredient(s): Pitavastatin calcium hydrate/Ezetimibe.
Notes: New combination drugs indicated for the treatment of hypercholesterolemia and familial hypercholesterolemia.